PMDA approval: Lumicef Subcutaneous Injection 210 mg Syringe
Applicant: Kyowa Kirin Co., Ltd. Approval date: 2020-11-27. Type: Partial change. Active ingredient(s): Brodalumab (genetical recombination).
Notes: A drug with new indications for the treatment of ankylosing spondylitis and non-radiographic axial spondyloarthritis in patients who have not responded sufficiently to conventional treatments.